PMDA approval: Xeloda Tablet 300
Applicant: Chugai Pharmaceutical Co., Ltd. Approval date: 2011-02-23. Type: Partial change. Active ingredient(s): Capecitabine.
Notes: A drug with a new additional indication and a new dosage for the treatment of unresectable advanced or recurrent gastric cancer. [Public knowledge-based application after PAFSC's preliminary assessment]